PMDA approval: Eylea 8 mg Solution for IVT Inj. 114.3 mg/mL
Applicant: Bayer Yakuhin, Ltd. Approval date: 2024-01-18. Type: New approval. Active ingredient(s): Aflibercept (genetical recombination).
Notes: A drug in a new dosage form indicated for the treatment of age-related macular degeneration associated with subfoveal choroidal neovascularization and diabetic macular edema.